PMDA approval: Alesion LX Ophthalmic Solution 0.1%
Applicant: Santen Pharmaceutical Co., Ltd. Approval date: 2019-09-20. Type: New approval. Active ingredient(s): Epinastine hydrochloride.
Notes: A drug with a new dosage in an additional dosage form indicated for the treatment of allergic conjunctivitis.